PMDA approval: Firdapse Tablets 10 mg
Applicant: DyDo Pharma, Inc. Approval date: 2024-09-24. Type: New approval. Active ingredient(s): Amifampridine phosphate.
Notes: A drug with a new active ingredient indicated for the improvement of muscle weakness in patients with Lambert-Eaton myasthenic syndrome. [Orphan drug]